PMDA approval: Inavir Dry Powder Inhaler 20 mg
Applicant: Daiichi Sankyo Company, Limited. Approval date: 2010-09-10. Type: New approval. Active ingredient(s): Laninamivir octanoate hydrate.
Notes: A drug with a new active ingredient indicated for the treatment of influenza A or B virus infections.